PMDA approval: Hympavzi S.C. Injection 150 mg Pen
Applicant: Pfizer Japan Inc. Approval date: 2024-12-27. Type: New approval. Active ingredient(s): Marstacimab (genetical recombination).
Notes: A drug with a new active ingredient indicated for the control of bleeding tendency in patients with congenital haemophilia who do not have inhibitors against blood coagulation factor VIII or IX.